PMDA approval: Tibsovo Tablets 250 mg
Applicant: Nihon Servier Co., Ltd. Approval date: 2025-03-27. Type: New approval. Active ingredient(s): Ivosidenib.
Notes: A drug with a new active ingredient indicated for the treatment of IDH1 mutation-positive acute myeloid leukemia. [Orphan drug]